PMDA approval: Treakisym Injection Solution 100 mg/4 mL
Applicant: SymBio Pharmaceuticals Limited. Approval date: 2022-02-25. Type: Partial change. Active ingredient(s): Bendamustine hydrochloride hydrate.
Notes: A drug with a new dosage indicated for the treatment of low-grade B-cell non-Hodgkin’s lymphoma, mantle cell lymphoma, chronic lymphocytic leukemia, and relapsed or refractory diffuse large B-cell lymphoma.